PMDA approval: Nexium Capsules 10 mg Nexium Capsules 20 mg Nexium Granules for Suspension 10 mg Nexium Granules for Suspension 20 mg
Applicant: AstraZeneca K.K. Approval date: 2024-06-24. Type: Partial change. Active ingredient(s): Esomeprazole magnesium hydrate.
Notes: Drugs with a new additional pediatric dosage indicated for the prevention of recurrent reflux esophagitis and gastric or duodenal ulcer after dosing non-steroid anti-inflammatory drugs, and prevention of recurrent gastric or duodenal ulcer after dosing low-dose aspirins.